PMDA approval: Adcetris for Intravenous Drip Infusion 50 mg
Applicant: Takeda Pharmaceutical Company Limited. Approval date: 2014-01-17. Type: New approval. Active ingredient(s): Brentuximab vedotin (genetical recombination).
Notes: A drug with a new active ingredient indicated for the treatment of relapsed or refractory CD30-positive Hodgkin’s lymphoma and anaplastic large-cell lymphoma. [Orphan drug]